PMDA approval: Selincro Tablets 10 mg
Applicant: Otsuka Pharmaceutical Co., Ltd. Approval date: 2019-01-08. Type: New approval. Active ingredient(s): Nalmefene hydrochloride hydrate.
Notes: A drug with a new active ingredient indicated to reduce alcohol consumption in patients with alcohol dependence.